PMDA approval: Scandonest Cartridge 3%
Applicant: Nippon Shika Yakuhin Co., Ltd. Approval date: 2022-12-23. Type: Partial change. Active ingredient(s): Mepivacaine hydrochloride.
Notes: A drug with a new indication for nerve block in the fields of dentistry and oral surgery. [Public knowledge-based application after PAFSC's preliminary assessment]